PMDA approval: Clarith Tab. 200 (Taisho Pharmaceutical Co., Ltd.) Klaricid Tablets 200 mg
Applicant: Abbott Japan Co., Ltd. Approval date: 2008-08-29. Type: Partial change. Active ingredient(s): Clarithromycin.
Notes: Drugs with a new indication and a new dosage for non-tuberculous mycobacteriosis including mycobacterium avium complex (MAC) infections.